PMDA approval: Imbruvica Capsules 140 mg
Applicant: Janssen Pharmaceutical K.K. Approval date: 2023-02-24. Type: Partial change. Active ingredient(s): Ibrutinib.
Notes: A drug with a new indication and a new dosage for the treatment of mantle cell lymphoma. [Orphan drug]